PMDA approval: Mirena 52 mg
Applicant: Bayer Yakuhin, Ltd. Approval date: 2014-06-20. Type: Partial change. Active ingredient(s): Levonorgestrel.
Notes: A drug with a new additional indication for the treatment of hypermenorrhea. [Public knowledge-based application after PAFSC's preliminary assessment]